PMDA approval: Fludara for IV Inj. 50 mg
Applicant: Bayer Yakuhin, Ltd. Approval date: 2009-11-06. Type: Partial change. Active ingredient(s): Fludarabine phosphate.
Notes: A drug with new additional indications and a new dosage for the treatment of recurrent or refractory low-grade B-cell non-Hodgkin’s lymphoma and mantle cell lymphoma.